PMDA approval: Rapalimus Tablets 1 mg
Applicant: Nobelpharma Co., Ltd. Approval date: 2021-09-27. Type: Partial change. Active ingredient(s): Sirolimus.
Notes: A drug with a new indication and a new dosage for the treatment of refractory lymphatic disease (lymphangioma [lymphatic malformation], lymphangiomatosis, Gorham's disease, and lymphangiectasia). [Orphan drug]